PMDA approval: Metreleptin for Subcutaneous Injection 11.25 mg "Shionogi"
Applicant: Shionogi & Co., Ltd. Approval date: 2013-03-25. Type: New approval. Active ingredient(s): Metreleptin (genetical recombination).
Notes: A drug with a new active ingredient for the treatment of lipodystrophy. [Orphan drug]